PMDA approval: Xofigo Injection
Applicant: Bayer Yakuhin, Ltd. Approval date: 2016-03-28. Type: New approval. Active ingredient(s): Radium （223Ra） dichloride.
Notes: A drug with a new active ingredient indicated for the treatment of castration-resistant prostate cancer with bone metastases.